PMDA approval: Viviant Tablets 20 mg
Applicant: Pfizer Japan Inc. Approval date: 2010-07-23. Type: New approval. Active ingredient(s): Bazedoxifene acetate.
Notes: A drug with a new active ingredient indicated for the treatment of postmenopausal osteoporosis.